PMDA approval: Botox for Injection 50 Units Botox for Injection 100 Units
Applicant: GlaxoSmithKline K.K. Approval date: 2019-12-20. Type: Partial change. Active ingredient(s): Botulinum toxin type A.
Notes: Drugs with a new route of administration indicated for the treatment of urinary urgency, urinary frequency, and urge urinary incontinence associated with overactive bladder or urinary incontinence due to neurogenic bladder, in patients who have not responded sufficiently to conventional therapies or in whom conventional therapies are not suitable.